PMDA approval: Strattera Capsules 5 mg Strattera Capsules 10 mg Strattera Capsules 25 mg
Applicant: Eli Lilly Japan K.K. Approval date: 2009-04-22. Type: New approval. Active ingredient(s): Atomoxetine hydrochloride.
Notes: Drugs with a new active ingredient indicated for the treatment of attention-deficit/hyperactivity disorder (AD/HD) in children. [Expedited review]